PMDA approval: Lunabell Tablets LD
Applicant: Nobelpharma Co., Ltd. Approval date: 2013-05-16. Type: Partial change. Active ingredient(s): Norethisterone/ ethinylestradiol.
Notes: A drug with a revised indication from "dysmenorrhea associated with endometriosis; functional dysmenorrhea" to "dysmenorrhea."